PMDA approval: Gla-alpha Combination Ophthalmic Solution
Applicant: Kowa Company, Ltd. Approval date: 2022-09-26. Type: New approval. Active ingredient(s): Ripasudil hydrochloride hydrate, Brimonidine tartrate.
Notes: A new combination drug indicated for the treatment of glaucoma and ocular hypertension in patients who have not responded sufficiently to other anti-glaucoma drugs.